PMDA approval: Mekinist Tablets 0.5 mg Mekinist Tablets 2 mg
Applicant: Novartis Pharma K.K. Approval date: 2016-03-28. Type: New approval. Active ingredient(s): Trametinib dimethyl sulfoxide.
Notes: Drugs with a new active ingredient indicated for the treatment of unresectable melanoma with BRAF mutation. [Orphan drug]